PMDA approval: NovoRapid 300 NovoRapid 300 FlexPen NovoRapid 100 U/mL Vial NovoRapid 30 Mix NovoRapid 30 Mix FlexPen
Applicant: Novo Nordisk Pharma Ltd. Approval date: 2007-08-23. Type: New approval. Active ingredient(s): Insulin aspart (genetical recombination).
Notes: Drugs containing a new active ingredient indicated for the treatment of diabetes mellitus where insulin therapy is indicated. A change has been made to the manufacturing process of the drug substance. Pharmaceutical formulations, manufacturing processes, indications, and dosage are the same as those in the approved formulations.